PMDA approval: Somatropin BS S.C. Injection 5 mg [Sandoz］ Somatropin BS S.C. Injection 10 mg [Sandoz]
Applicant: Sandoz K.K. Approval date: 2009-06-22. Type: New approval. Active ingredient(s): Somatropin (genetical recombination).
Notes: Follow-on biologics indicated for the treatment of growth disturbance due to growth hormone deficiency before epiphyseal closure and growth disturbance associated with Turner syndrome or chronic renal insufficiency before epiphyseal closure.